PMDA approval: Vtama Cream 1%
Applicant: Japan Tobacco Inc. Approval date: 2024-06-24. Type: New approval. Active ingredient(s): Tapinarof.
Notes: A drug with a new active ingredient indicated for the treatment of atopic dermatitis and plaque psoriasis.